PMDA approval: mRESVIA Intramuscular Injection Syringes
Applicant: Moderna Japan Co., Ltd. Approval date: 2025-05-19. Type: New approval. Active ingredient(s): mRNA encoding the F glycoprotein from RSV subtype A stabilized in the prefusion conformation.
Notes: A drug with a new active ingredient indicated for the prevention of disease caused by RS virus infection.